PMDA approval: Opdivo Intravenous Infusion 20 mg Opdivo Intravenous Infusion 100 mg
Applicant: Ono Pharmaceutical Co., Ltd. Approval date: 2017-03-24. Type: Partial change. Active ingredient(s): Nivolumab (genetical recombination).
Notes: Drugs with a new additional indication for the treatment of relapse or distant metastasis of head and neck cancer. [Priority review]